PMDA approval: Xolair for S.C. Injection 150 mg Xolair for S.C. Injection 75 mg
Applicant: Novartis Pharma K.K. Approval date: 2013-08-20. Type: Partial change. Active ingredient(s): Omalizumab (genetical recombination).
Notes: Drugs with a new dosage indicated for the treatment of bronchial asthma (for use only in patients with intractable bronchial asthma whose asthmatic responses are uncontrollable with conventional therapies).